PMDA approval: Retympa 250 μg Set for Otology
Applicant: Nobelpharma Co., Ltd. Approval date: 2019-09-20. Type: New approval. Active ingredient(s): Trafermin (genetical recombination).
Notes: A drug with a new route of administration indicated for the treatment of tympanic membrane perforation.